PMDA approval: Divigel 1 mg
Applicant: Sun Pharma Japan Ltd. Approval date: 2022-03-11. Type: Partial change. Active ingredient(s): Estradiol.
Notes: A drug with a new indication and a new dosage for adjusting the start timing of controlled ovarian stimulation in assisted reproductive technology and hormone replacement cycle in frozen-thawed embryo transfer. [Public knowledge-based application]